PMDA approval: Tauvid Injection
Applicant: PDRadiopharma Inc. Approval date: 2024-12-27. Type: New approval. Active ingredient(s): Flortaucipir (18F).
Notes: A drug with a new active ingredient indicated for the support of selection of patients with mild cognitive impairment and mild dementia due to Alzheimer's disease, for whom donanemab (genetical recombination) is indicated. [Expedited review]